PMDA approval: Nexavar Tablets 200 mg
Applicant: Bayer Yakuhin, Ltd. Approval date: 2009-05-20. Type: Partial change. Active ingredient(s): Sorafenib tosilate.
Notes: A drug with a new additional indication for the treatment of unresectable hepatocellular carcinoma. [Priority review]